PMDA approval: Marduox Ointment
Applicant: Chugai Pharmaceutical Co., Ltd. Approval date: 2016-03-28. Type: New approval. Active ingredient(s): Maxacalcitol / Betamethasone butyrate propionate.
Notes: A new combination drug indicated for the treatment of psoriasis vulgaris.